PMDA approval: Vyvgart for Intravenous Infusion 400 mg
Applicant: Argenx Japan K.K. Approval date: 2022-01-20. Type: New approval. Active ingredient(s): Efgartigimod alfa (genetical recombination).
Notes: A drug with a new active ingredient indicated for the treatment of generalized myasthenia gravis (for use only in patients who have not sufficiently responded to steroids or other immunosuppressants). [Orphan drug]